PMDA approval: Dormicum Syrup 2 mg/mL
Applicant: Maruishi Pharmaceutical Co., Ltd. Approval date: 2025-09-19. Type: New approval. Active ingredient(s): Midazolam.
Notes: A drug with a new route of administration indicated for anesthetic premedication. [Drugs for specific use]